PMDA approval: Remeron Tablets 15 mg (Schering-Plough K.K.) Reflex Tablets 15 mg
Applicant: Meiji Seika Kaisha, Ltd. Approval date: 2009-07-07. Type: New approval. Active ingredient(s): Mirtazapine.
Notes: Drugs with a new active ingredient indicated for the treatment of depression.